PMDA approval: Apohide Lotion 20%
Applicant: Hisamitsu Pharmaceutical Co., Inc. Approval date: 2023-03-27. Type: New approval. Active ingredient(s): Oxybutynin hydrochloride.
Notes: A drug with a new indication in a new dosage form for the treatment of primary palmar hyperhidrosis.